PMDA approval: Dupixent 300 mg Syringe for S.C. Injection
Applicant: Sanofi K.K. Approval date: 2020-03-25. Type: Partial change. Active ingredient(s): Dupilumab (genetical recombination).
Notes: A drug with a new additional indication and a new dosage for the treatment of chronic rhinosinusitis with nasal polyps (for use only in patients who have not responded sufficiently to conventional treatments).